PMDA approval: Emulsion-adjuvanted Cell-culture Derived Influenza HA Vaccine H5N1 for Intramuscular Injection “Kaketsuken”
Applicant: Kaketsuken [The Chemo-Sero-Therapeutic Research Institute]. Approval date: 2018-03-23. Type: Partial change. Active ingredient(s): Emulsion-adjuvanted cell-culture derived influenza HA vaccine (H5N1).
Notes: A drug with a new additional pediatric dosage indicated for the prevention of pandemic influenza (H5N1). [Orphan drug]